PMDA approval: Miripla Suspension Vehicle 4 mL
Applicant: Dainippon Sumitomo Pharma Co., Ltd. Approval date: 2009-08-20. Type: New approval. Active ingredient(s): Iodine addition products of the ethyl esters of the fatty acids obtained from poppyseed oil.
Notes: A drug with a new indication for suspending Miripla for intra-arterial injection 70 mg.